PMDA approval: Onbrez Inhalation Capsules 150 μg
Applicant: Novartis Pharma K.K. Approval date: 2011-07-01. Type: New approval. Active ingredient(s): Indacaterol maleate.
Notes: A drug with a new active ingredient indicated for the alleviation of various symptoms due to airway obstructive impairment in chronic obstructive pulmonary diseases (chronic bronchitis and emphysema).